PMDA approval: Galafold Capsules 123 mg
Applicant: Amicus Therapeutics, Inc. Approval date: 2018-03-23. Type: New approval. Active ingredient(s): Migalastat hydrochloride.
Notes: A drug with a new active ingredient indicated for the treatment of Fabry disease in patients with GLA mutations categorized as amenable to treatment with migalastat. [Orphan drug]